PMDA approval: Xiaflex lnj.
Applicant: Asahi Kasei Pharma Corporation. Approval date: 2015-07-03. Type: New approval. Active ingredient(s): Collagenase (Clostridium histolyticum ).
Notes: A drug with a new active ingredient indicated for the treatment of Dupuytren's contracture.